PMDA approval: Tivicay Tablets 50 mg
Applicant: ViiV Healthcare K.K. Approval date: 2014-03-24. Type: New approval. Active ingredient(s): Dolutegravir sodium.
Notes: A drug with a new active ingredient indicated for the treatment of HIV infection. [Orphan drug]